PMDA approval: Ilumya Subcutaneous Injection 100 mg Syringe
Applicant: Sun Pharma Japan Ltd. Approval date: 2020-06-29. Type: New approval. Active ingredient(s): Tildrakizumab (genetical recombination).
Notes: A drug with a new active ingredient indicated for the treatment of plaque psoriasis in patients who have not responded sufficiently to conventional therapies.